PMDA approval: Revlimid Capsules 5 mg
Applicant: Celgene K.K. Approval date: 2010-08-20. Type: Partial change. Active ingredient(s): Lenalidomide hydrate.
Notes: A drug with a new additional indication and a new dosage for the treatment of myelodysplastic syndrome associated with a deletion 5q cytogenetic abnormality. [Orphan drug]